PMDA approval: Sovaldi Tablets 400 mg
Applicant: Gilead Sciences, Inc. Approval date: 2017-03-24. Type: Partial change. Active ingredient(s): Sofosbuvir.
Notes: A drug with a new additional indication and a new dosage for the improvement of viraemia in patients with chronic hepatitis C or compensated cirrhosis type C in neither Serogroup 1 (genotype 1) nor Serogroup 2 (genotype 2). [Expedited review]